PMDA approval: Lyrica Capsules 25 mg Lyrica Capsules 75 mg Lyrica Capsules 150 mg
Applicant: Pfizer Japan Inc. Approval date: 2012-06-22. Type: Partial change. Active ingredient(s): Pregabalin.
Notes: Drugs with a new additional indication and a new dosage for the treatment of pain associated with fibromyalgia. [Priority review]